PMDA approval: Taxol Injection 30 mg Taxol Injection 100 mg (Bristol-Myers K.K.) Paclitaxel Inj. 30 mg/5 mL "NK" Paclitaxel Inj. 100 mg/16.7 mL "NK" (Nippon Kayaku Co., Ltd.) Paclitaxel I.V. Infusion 30 mg/5 mL "Hospira" Paclitaxel I.V. Infusion 100 mg/16.7 mL "Hospira" (Hospira Japan Co., Ltd.) Paclitaxel Injection 30 mg "Sawai" Paclitaxel Injection 100 mg "Sawai" Paclitaxel Injection 150 mg "Sawai" (Sawai Pharmaceutical Co., Ltd.) Paclitaxel Intravenous Infusion 30 mg "Sandoz" Paclitaxel Intravenous Infusion 100 mg "Sandoz" (Sandoz K.K.) Paclitaxel Injection 30 mg "NP" Paclitaxel Injection 100 mg "NP" (Nipro Corporation) Paclitaxel Injection 30 mg "Pfizer" Paclitaxel Injection 100 mg "Pfizer"
Applicant: Mylan Seiyaku Ltd. Approval date: 2015-09-24. Type: Partial change. Active ingredient(s): Paclitaxel.
Notes: Drugs with a new dosage indicated for the treatment of gastric cancer. [Public knowledge-based application after PAFSC's preliminary assessment]